PMDA approval: Encevac for Subcutaneous Injection
Applicant: Kaketsuken [The Chemo-Sero-Therapeutic Research Institute]. Approval date: 2011-01-17. Type: New approval. Active ingredient(s): Freeze-dried, cell culture-derived Japanese encephalitis vaccine （inactivated）.
Notes: A drug with a new active ingredient indicated for the prevention of Japanese encephalitis. [Expedited review]